PMDA approval: MabCampath Intravenous infusion 30 mg
Applicant: Sanofi K.K. Approval date: 2014-09-26. Type: New approval. Active ingredient(s): Alemtuzumab (genetical recombination).
Notes: A drug with a new active ingredient indicated for the treatment of relapsed or refractory chronic lymphocytic leukemia. [Orphan drug]